PMDA approval: Fentos Tape 0.5 mg Fentos Tape 1 mg Fentos Tape 2 mg Fentos Tape 4 mg Fentos Tape 6 mg Fentos Tape 8 mg
Applicant: Hisamitsu Pharmaceutical Co., Inc. Approval date: 2021-08-25. Type: Partial change. Active ingredient(s): Fentanyl citrate.
Notes: Drugs with a new indication and a new additional pediatric dosage for the analgesia in various types of cancer with moderate to severe pain that cannot be managed with non-opioid analgesics (for use only in patients who switch from other opioid analgesics).